PMDA approval: Shingrix for I.M. Injection
Applicant: GlaxoSmithKline K.K. Approval date: 2023-06-26. Type: Partial change. Active ingredient(s): Varicella zoster virus gE antigen.
Notes: A drug with a new dosage indicated for the prevention of herpes zoster.